PMDA approval: Duac Combination Gel
Applicant: GlaxoSmithKline K.K. Approval date: 2015-03-26. Type: New approval. Active ingredient(s): Clindamycin phosphate hydrate/ Benzoyl peroxide.
Notes: A new combination drug with a new active ingredient indicated for the treatment of acne vulgaris.